PMDA approval: Rapifort Wipes 2.5%
Applicant: Maruho Co., Ltd. Approval date: 2022-01-20. Type: New approval. Active ingredient(s): Glycopyrronium tosilate hydrate.
Notes: A drug with a new active ingredient indicated for the treatment of primary axillary hyperhidrosis.